PMDA approval: Entresto Tablets 100 mg Entresto Tablets 200 mg
Applicant: Novartis Pharma K.K. Approval date: 2021-09-27. Type: Partial change. Active ingredient(s): Sacubitril valsartan sodium hydrate.
Notes: Drugs with a new indication and a new dosage for the treatment of hypertension.